PMDA approval: Juluca Combination Tablets
Applicant: ViiV Healthcare K.K. Approval date: 2018-11-26. Type: New approval. Active ingredient(s): Dolutegravir sodium/Rilpivirine hydrochloride.
Notes: A new combination drug with a new active ingredient indicated for the treatment of HIV-1 infection. [Orphan drug]